PMDA approval: Mycobutin Capsules 150 mg
Applicant: Pfizer Japan Inc. Approval date: 2008-07-16. Type: New approval. Active ingredient(s): Rifabutin.
Notes: A drug containing a new active ingredient indicated for the treatment of 1) tuberculosis, 2) non-tuberculous mycobacteriosis including mycobacterium avium complex (MAC) infections, and 3) prophylaxis of disseminated MAC infections in patients with HIV disease.